PMDA approval: Repatha･SC Injection 140 mg Syringe Repatha･SC Injection 140 mg Pen
Applicant: Amgen Astellas BioPharma K.K. Approval date: 2016-01-22. Type: New approval. Active ingredient(s): Evolocumab (genetical recombination).
Notes: Drugs with a new active ingredient indicated for the treatment of familial hypercholesterolemia and hypercholesterolemia (for use only in patients who are at higher risk of developing cardiovascular event and have not responded sufficiently to HMG-CoA reductase inhibitors).